PMDA approval: Xalkori Capsules 200 mg Xalkori Capsules 250 mg
Applicant: Pfizer Japan Inc. Approval date: 2012-03-30. Type: New approval. Active ingredient(s): Crizotinib.
Notes: Drugs with a new active ingredient indicated for the treatment of unresectable advanced/relapsed ALK fusion gene-positive non-small-cell lung cancer. [Orphan drug]